PMDA approval: Darbepoetin Alfa BS Injection 5 μg Syringe [JCR] Darbepoetin Alfa BS Injection 10 μg Syringe [JCR] Darbepoetin Alfa BS Injection 15 μg Syringe [JCR] Darbepoetin Alfa BS Injection 20 μg Syringe [JCR] Darbepoetin Alfa BS Injection 30 μg Syringe [JCR] Darbepoetin Alfa BS Injection 40 μg Syringe [JCR] Darbepoetin Alfa BS Injection 60 μg Syringe [JCR] Darbepoetin Alfa BS Injection 120 μg Syringe [JCR] Darbepoetin Alfa BS Injection 180 μg Syringe [JCR]
Applicant: JCR Pharmaceuticals Co., Ltd. Approval date: 2019-09-20. Type: New approval. Active ingredient(s): Darbepoetin alfa (genetical recombination) [darbepoetin alfa biosimilar 1].
Notes: Follow-on biologics indicated for the treatment of nephrogenic anaemia.